PMDA approval: Prograf Capsules 0.5 mg Prograf Capsules 1 mg Prograf Granules 0.2 mg Prograf Granules 1 mg
Applicant: Astellas Pharma Inc. Approval date: 2009-10-16. Type: Partial change. Active ingredient(s): Tacrolimus hydrate.
Notes: Drugs with a revised indication for the treatment of myasthenia gravis (limitation of patients to be treated was abolished.) [Orphan drug]